PMDA approval: Comirnaty Intramuscular Injection for 5 to 11 years old
Applicant: Pfizer Japan Inc. Approval date: 2023-02-28. Type: Partial change. Active ingredient(s): Tozinameran Tozinameran and Famtozinameran.
Notes: A drug with a new indication, a new dosage, and other characteristics for the prevention of disease caused by SARS-CoV-2 infection (COVID-19). [Drug pertaining to Special Approval for Emergency]